PMDA approval: Singulair Fine Granules 4 mg (Banyu Pharmaceutical Co., Ltd.) Kipres Fine Granules 4 mg
Applicant: Kyorin Pharmaceutical Co., Ltd. Approval date: 2007-07-31. Type: New approval. Active ingredient(s): Montelukast sodium.
Notes: Drugs with a new dosage and in a new dosage form, or fine granules, indicated for the treatment of asthma in children aged 1 or more and less than 6 years. 6-1 53 Calonal Fine Granules 20% Change Acetaminophen Calonal Fine Granules 50% Change Calonal Tablets 200 Change Calonal Tablets 300 Change Calonal Syrup 2% Change Calonal Suppositories 100 Change Calonal Suppositories 200 Change (Showa Yakuhin Kako Co., Ltd.) Pyrinazin Powder Change (Astellas Pharma Inc.) Anhiba Suppositories 50 mg for Pediatric Use Change Anhiba Suppositories 100 mg for Pediatric Use Change Anhiba Suppositories 200 mg for Pediatric Use Change (Abbott Japan Co., Ltd.) Pyretinol Change (Iwaki Seiyaku Co.,Ltd.) Acetaminophen Tablets 200 mg "Isei" Change (Isei Co., Inc.) Cocarl Dry Syrup 20% for Pediatric Change Cocarl Dry Syrup 40% Change Cocarl Tablets 200 mg Change (Sanwa Kagaku Kenkyusho Co., Ltd.) Paraceta Suppositories 100 Change Paraceta Suppositories 200 Change (Sioe Pharmaceutical Co., Ltd.) Acetaminophen Tablets 200 mg (TYK) Change Acetaminophen Suppositories 100 Change Acetaminophen Suppositories 200 Change Acetaminophen Fine Granules 20% (TYK) Change (Taisho Pharmaceutical Co., Ltd.) Calsil Fine Granules 20% Change Calsil Tablets 200 Change Calsil Syrup 2% for Pediatric Use Change Calsil Suppositories 200 for Pediatric Use Change Sep. 28, 2007 Drugs with a new indication and dosage for use as an antipyretic/analgesic in pediatric patients. Expedited review. Calsil Suppositories 100 for Pediatric Use Change (Taiyo Yakuhin Co., Ltd.) Atmiphen Tablets 200 Change Atmiphen Dry Syrup 20% Change (Takata Seiyaku Co., Ltd.) Acetaminophen Tablets 200 "Tatsumi" Change Acetaminophen Fine Granules 20% "Tatsumi" Change (Tatsumi Kagaku Co., Ltd.) Anyrume Tablets 200 mg Change Anyrume Tablets 300 mg Change Anyrume S Change Anyrume S Suppositories 200 Change Anyrume Fine Granules Change (Choseido Pharmaceutical Co., Ltd.) Atenemen 20% Fine Granules Change Atenemen Tablets 200 mg Change Atenemen Suppositories 100 mg Change Atenemen Suppositories 200 mg Change (Tsuruhara Pharmaceutical Co., Ltd.) Acetaminophen "Hachi" Change (Oriental Pharmaceutical and Synthetic Chemical Co., Ltd.) Sarutu Fine Granules 20% Change Sarutu Tablets 200 mg Change Sarutu Syrup for Pediatric 2% Change Sarutu Dry Syrup for Pediatric 20% Change (Towa Pharmaceutical Co., Ltd.) Aphlogis Suppositories 100 Change Aphlogis Suppositories 200 Change (Nissin Pharmaceutical Co., Ltd. ) Acetaminophen Tablets 200 mg "NP" Change Acetaminophen Fine Granules 20% Change (Nipro Pharma Corporation) Alpiny Suppositories 50 Change Alpiny Suppositories 100 Change Alpiny Suppositories 200 Change (Hisamitsu Pharmaceutical Co., Inc.) Aspain Change (Maruishi Pharmaceutical Co., Ltd.) Napa Change Napa Dry Syrup 20% Change (Merck Seiyaku Ltd.) Acetaminophen "Yoshida" Change (Yoshida Pharmaceutical Co., Ltd.)